PMDA approval: Venoglobulin IH 10% I.V. 0.5 g/5 mL Venoglobulin IH 10% I.V. 2.5 g/25 mL Venoglobulin IH 10% I.V. 5 g/50 mL Venoglobulin IH 10% I.V. 10 g/100 mL Venoglobulin IH 10% I.V. 20 g/200 mL
Applicant: Japan Blood Products Organization. Approval date: 2024-09-24. Type: Partial change. Active ingredient(s): Polyethylene glycol treated human normal immunoglobulin.
Notes: Drugs with new indications and a new dosage for the treatment of antibody-mediated rejection in the following organ transplantations; kidney transplantation, liver transplantation, heart transplantation, lung transplantation, pancreas transplantation, small intestine transplantation. [Orphan drug]